PMDA approval: Hycamtin for Injection 1.1 mg
Applicant: Nippon Kayaku Co., Ltd. Approval date: 2013-06-14. Type: Partial change. Active ingredient(s): Nogitecan hydrochloride.
Notes: A drug with a new additional indication and a new dosage for the treatment of pediatric malignant solid tumors. [Public knowledge-based application after PAFSC's preliminary assessment]